PMDA approval: Enbrel 25 mg for S.C. Injection Enbrel 25 mg Syringe 0.5 mL for S.C. Injection Enbrel 10 mg for S.C. Injection Enbrel 50 mg Syringe 1.0 mL for S.C. Injection
Applicant: Pfizer Japan Inc. Approval date: 2012-03-21. Type: Partial change. Active ingredient(s): Etanercept (genetical recombination).
Notes: Drugs with a new additional indication for the treatment of rheumatoid arthritis (including prevention of structural joint damage) in patients who have not sufficiently responded to conventional treatments.